PMDA approval: Nasonex Nasal Suspension 50 μg 56 spray Nasonex Nasal Suspension 50 μg 112 spray
Applicant: Schering-Plough K.K. Approval date: 2008-07-16. Type: New approval. Active ingredient(s): Mometasone furoate hydrate.
Notes: Drugs containing a new active ingredient indicated for the treatment of allergic rhinitis.